PMDA approval: Orencia SC 125 mg Syringe 1 mL
Applicant: Bristol-Myers K.K. Approval date: 2013-06-28. Type: New approval. Active ingredient(s): Abatacept (genetical recombination).
Notes: A drug with a new route of administration and a new dosage in a new dosage form indicated for the treatment of rheumatoid arthritis in patients who have not responded sufficiently to conventional treatments.